PMDA approval: Rosuzet Combination Tablets LD Rosuzet Combination Tablets HD
Applicant: MSD K.K. Approval date: 2019-03-26. Type: New approval. Active ingredient(s): Ezetimibe/Rosuvastatin calcium.
Notes: New combination drugs indicated for the treatment of hypercholesterolemia and familial hypercholesterolemia.